PMDA approval: Venoglobulin IH 5% I.V. 2.5 g/50 mL
Applicant: Japan Blood Products Organization. Approval date: 2015-02-02. Type: Partial change. Active ingredient(s): Polyethylene glycol treated human normal immunoglobulin.
Notes: A drug with a new indication and a new dosage for the prevention of acute otitis media, acute bronchitis, or pneumonia caused by Pneumococcus or Haemophilus influenzae in patients associated with a decrease in serum IgG2 levels. (for use only in patients who have not responded sufficiently to prevention by vaccination and other appropriate treatments, and have repeated the relapse of these diseases).